PMDA approval: Fludara 50 mg for Intravenous Infusion
Applicant: Sanofi K.K. Approval date: 2019-03-26. Type: Partial change. Active ingredient(s): Fludarabine phosphate.
Notes: A drug with other characteristics indicated for the treatment prior to tumor-specific T-cell infusion therapy. [Expedited review]